PMDA approval: Recalbon Tablets 1 mg (Ono Pharmaceutical Co., Ltd.) Bonoteo Tablets 1 mg
Applicant: Astellas Pharma Inc. Approval date: 2009-01-21. Type: New approval. Active ingredient(s): Minodronic acid hydrate.
Notes: Drugs containing a new active ingredient indicated for the treatment of osteoporosis.